PMDA approval: Piasky for Injection 340 mg
Applicant: Chugai Pharmaceutical Co., Ltd. Approval date: 2024-03-26. Type: New approval. Active ingredient(s): Crovalimab (genetical recombination).
Notes: A drug with a new active ingredient indicated for the treatment of paroxysmal nocturnal haemoglobinuria.